PMDA approval: Smyraf Tablets 50 mg Smyraf Tablets 100 mg
Applicant: Astellas Pharma Inc. Approval date: 2019-03-26. Type: New approval. Active ingredient(s): Peficitinib hydrobromide.
Notes: Drugs with a new active ingredient indicated for the treatment of rheumatoid arthritis (including prevention of structural joint damage) in patients who have not responded sufficiently to conventional treatments.